PMDA approval: Unitalc Intrapleural 4 g
Applicant: Nobelpharma Co., Ltd. Approval date: 2022-03-28. Type: Partial change. Active ingredient(s): Talc.
Notes: A drug with a new indication for the treatment of inoperable secondary intractable pneumothorax. [Orphan drug]